PMDA approval: E Keppra Tablets 250 mg E Keppra Tablets 500 mg
Applicant: UCB Japan Co., Ltd. Approval date: 2013-05-31. Type: Partial change. Active ingredient(s): Levetiracetam.
Notes: Drugs with a new additional pediatric dosage indicated for use as an adjunctive therapy with other antiepileptic drugs to treat partial seizures (including secondary generalized seizures) in patients with epilepsy who have not responded sufficiently to other antiepileptic drugs.